PMDA approval: Bavencio Injection 200 mg
Applicant: Merck Serono Co., Ltd. Approval date: 2017-09-27. Type: New approval. Active ingredient(s): Avelumab (genetical recombination).
Notes: A drug with a new active ingredient indicated for the treatment of unresectable Merkel cell carcinoma. [Orphan drug]